PMDA approval: Tramal Capsules 25 mg Tramal Capsules 50 mg
Applicant: Nippon Shinyaku Co., Ltd. Approval date: 2013-06-14. Type: Partial change. Active ingredient(s): Tramadol hydrochloride.
Notes: Drugs with a new additional indication for analgesia in patients with chronic non-cancer pain which cannot be managed by treatments with non-opioid analgesics.